PMDA approval: Avastin 100 mg/4 mL Intravenous Infusion Avastin 400 mg/16 mL Intravenous Infusion
Applicant: Chugai Pharmaceutical Co., Ltd. Approval date: 2016-05-23. Type: Partial change. Active ingredient(s): Bevacizumab (genetical recombination).
Notes: Drugs with a new additional indication for the treatment of advanced or recurrent cervical cancer. [Orphan drug]